PMDA approval: Kenketu Nonthron 500 for injection Kenketu Nonthron 1500 for injection
Applicant: Nihon Pharmaceutical Co., Ltd. Approval date: 2017-08-25. Type: Partial change. Active ingredient(s): Lyophilized human antithrombin III concentrate.
Notes: Drugs with a new additional indication and a new dosage for the treatment of portal vein thrombosis associated with decreased antithrombin III.